PMDA approval: Menoaid Combipatch
Applicant: ASKA Pharmaceutical. Co., Ltd. Approval date: 2008-10-16. Type: New approval. Active ingredient(s): Estradiol/norethisterone acetate.
Notes: A new combination drug indicated for the treatment of vasomotor symptoms (hot flushes and sweating) associated with climacteric disturbance and ovarian deficiency symptoms.